PMDA approval: Remicade for I.V. Infusion 100
Applicant: Mitsubishi Tanabe Pharma Corporation. Approval date: 2010-04-16. Type: Partial change. Active ingredient(s): Infliximab (genetical recombination).
Notes: A drug with a new additional indication and a new dosage for the treatment of ankylosing spondylitis in patients who have not responded sufficiently to conventional treatments. [Orphan drug]